PMDA approval: Diquas Ophthalmic Solution 3%
Applicant: Santen Pharmaceutical Co., Ltd. Approval date: 2010-04-16. Type: New approval. Active ingredient(s): DiquafosoI sodium.
Notes: A drug with a new active ingredient indicated for the treatment of dry eye.